PMDA approval: Zepbound Subcutaneous Injection 2.5 mg Ateos Zepbound Subcutaneous Injection 5 mg Ateos Zepbound Subcutaneous Injection 7.5 mg Ateos Zepbound Subcutaneous Injection 10 mg Ateos Zepbound Subcutaneous Injection 12.5 mg Ateos Zepbound Subcutaneous Injection 15 mg Ateos
Applicant: Eli Lilly Japan K.K. Approval date: 2024-12-27. Type: New approval. Active ingredient(s): Tirzepatide.
Notes: Drugs with a new indication, a new dosage, and other characteristics for the treatment of obesity. For use only in patients with any of hypertension, hyperlipidemia or type 2 diabetes mellitus who have not responded sufficiently to diet therapy and exercise therapy, and meet the following conditions: • BMI of 27 kg/m2 or greater in the presence of at least two obesity-related comorbidities or • BMI of 35 kg/m2 or greater